PMDA approval: Popscaine 0.75% Inj. 75 mg/10 mL Popscaine 0.75% Inj. 150 mg/20 mL Popscaine 0.25% Inj. 25 mg/10 mL Popscaine 0.25% Inj. Bag 250 mg/100 mL Popscaine 0.75% Inj. Syringe 75 mg/10 mL Popscaine 0.25% Inj. Syringe 25 mg/10 mL
Applicant: Maruishi Pharmaceutical Co., Ltd. Approval date: 2008-04-16. Type: New approval. Active ingredient(s): Levobupivacaine hydrochloride.
Notes: Drugs containing a new active ingredient. The 0.25% formulation is indicated for postoperative analgesia, and the 0.75% formulation is indicated for epidural anaesthesia.